PMDA approval: (1) Wegovy Subcutaneous Injection 0.25 mg SD (1) Wegovy Subcutaneous Injection 0.5 mg SD (1) Wegovy Subcutaneous Injection 1.0 mg SD (2) Wegovy Subcutaneous Injection 1.7 mg SD (2) Wegovy Subcutaneous Injection 2.4 mg SD
Applicant: Novo Nordisk Pharma Ltd. Approval date: 2023-03-27. Type: New approval. Active ingredient(s): Semaglutide (genetical recombination).
Notes: (1) Drugs with a new indication, a new dosage and other characteristics for the treatment of obesity. (2) Drugs with a new indication and a new dosage in an additional dosage form for the treatment of obesity. For use only in patients with any of hypertension, hyperlipidemia or type 2 diabetes mellitus who have not responded sufficiently to diet therapy and exercise therapy, and meet the following conditions: • BMI of 27 kg/m2 or greater in the presence of at least two obesity-related comorbidities or • BMI of 35 kg/m2 or greater